PMDA approval: Vizamyl Injection
Applicant: Nihon Medi-Physics Co., Ltd. Approval date: 2023-08-31. Type: Partial change. Active ingredient(s): Flutemetamol (18F).
Notes: A drug with a new indication for the visualization of beta‑amyloid plaques in the brain of patients suspected of mild cognitive impairment or dementia due to Alzheimer's disease. [Expedited review]